PMDA approval: Opdivo Intravenous Infusion 20 mg Opdivo Intravenous Infusion 100 mg Opdivo Intravenous Infusion 120 mg Opdivo Intravenous Infusion 240 mg
Applicant: Ono Pharmaceutical Co., Ltd. Approval date: 2025-08-25. Type: Partial change. Active ingredient(s): Nivolumab (genetical recombination).
Notes: Drugs with a new indication and new dosage for the treatment of unresectable, advanced or recurrent microsatellite instability-high (MSI-High) colorectal cancer. [Orphan drug]